PMDA approval: Lunsumio for Intravenous Infusion 1 mg Lunsumio for Intravenous Infusion 30 mg
Applicant: Chugai Pharmaceutical Co., Ltd. Approval date: 2024-12-27. Type: New approval. Active ingredient(s): Mosunetuzumab (genetical recombination).
Notes: Drugs with a new active ingredient indicated for the treatment of relapsed or refractory follicular lymphoma.